PMDA approval: Zelboraf Tablet 240 mg
Applicant: Chugai Pharmaceutical Co., Ltd. Approval date: 2014-12-26. Type: New approval. Active ingredient(s): Vemurafenib.
Notes: A drug with a new active ingredient indicated for the treatment of unresectable malignant melanoma with BRAF mutation. [Orphan drug]